PMDA approval: Octreoscan Injection
Applicant: Fujifilm RI Pharma Co.,Ltd. Approval date: 2015-09-28. Type: New approval. Active ingredient(s): Indium pentetoreotide (111In).
Notes: A drug with a new active ingredient indicated for the diagnosis of neuroendocrine tumor by using somatostatin receptor scintigraphy.